PMDA approval: Decadron Tablets 0.5 mg Decadron Tablets 4 mg
Applicant: Nichi-Iko Pharmaceutical Co., Ltd. Approval date: 2021-08-25. Type: Partial change. Active ingredient(s): Dexamethasone.
Notes: Drugs with a new indication and a new dosage for the treatment of systemic AL amyloidosis. [Expedited review]